PMDA approval: Normosang Infusion 250 mg
Applicant: CMIC Holdings Co., Ltd. Approval date: 2013-03-25. Type: New approval. Active ingredient(s): Hemin.
Notes: A drug with a new active ingredient indicated for the improvement of symptoms of acute attack in patients with acute porphyria. [Orphan drug]